PMDA approval: Ajovy Syringes for S.C. Injection 225 mg
Applicant: Otsuka Pharmaceutical Co., Ltd. Approval date: 2021-06-23. Type: New approval. Active ingredient(s): Fremanezumab (genetical recombination).
Notes: A drug with a new active ingredient indicated for the prevention of migraine attacks.